PMDA approval: Ultomiris for intravenous Infusion 300 mg
Applicant: Alexion Pharma G.K. Approval date: 2019-06-18. Type: New approval. Active ingredient(s): Ravulizumab (genetical recombination).
Notes: A drug with a new active ingredient indicated for the treatment of paroxysmal nocturnal hemoglobinuria. [Orphan drug]